PMDA approval: Prevenar 13 Suspension Liquid for Injection
Applicant: Pfizer Japan Inc. Approval date: 2014-06-20. Type: Partial change. Active ingredient(s): Pneumococcal 13-valent conjugate vaccine adsorbed (mutated diphtheria CRM197 conjugate).
Notes: A drug with a new additional indication and a new dosage for the prophylaxis of pneumococcal disease. (serotypes 1, 3, 4, 5, 6A, 6B, 7F, 9V, 14, 18C, 19A, 19F, and 23F) in the elderly.